PMDA approval: Benlysta for I.V. Infusion 120 mg Benlysta for I.V. Infusion 400 mg
Applicant: GlaxoSmithKline K.K. Approval date: 2019-09-20. Type: Partial change. Active ingredient(s): Belimumab (genetical recombination).
Notes: Drugs with a new additional pediatric dosage indicated for the treatment of systemic lupus erythematosus in patients who have not responded sufficiently to conventional treatments.